PMDA approval: Haruropi Tape 8 mg Haruropi Tape 16 mg Haruropi Tape 24 mg Haruropi Tape 32 mg Haruropi Tape 40 mg
Applicant: Hisamitsu Pharmaceutical Co., Inc. Approval date: 2019-09-20. Type: New approval. Active ingredient(s): Ropinirole hydrochloride.
Notes: Drugs with a new route of administration indicated for the treatment of Parkinson's disease.